PMDA approval: Copaxone S.C. Injection 20 mg Syringe
Applicant: Takeda Pharmaceutical Company Limited. Approval date: 2015-09-28. Type: New approval. Active ingredient(s): Glatiramer acetate.
Notes: A drug with a new active ingredient indicated for the prevention of relapse in multiple sclerosis. [Orphan drug]